PMDA approval: Lonsurf Combination Tablet T15 Lonsurf Combination Tablet T20
Applicant: Taiho Pharmaceutical Co., Ltd. Approval date: 2019-08-22. Type: Partial change. Active ingredient(s): Trifluridine/Tipiracil hydrochloride.
Notes: Drugs with a new indication for the treatment of unresectable advanced or recurrent gastric cancer that have progressed after chemotherapy.